PMDA approval: Prevenar Suspension Liquid for S.C. Injection
Applicant: Wyeth K.K. Approval date: 2009-10-16. Type: New approval. Active ingredient(s): Pneumococcal polysaccharide (serotypes 4, 6B, 9V, 14, 18C, 19F, and 23F)-CRM197 conjugate.
Notes: A drug with a new active ingredient indicated for prophylaxis of pneumococcal invasive disease (serotypes 4, 6B, 9V, 14, 18C, 19F, and 23F). [Priority review]